PMDA approval: Keytruda Injection 100 mg
Applicant: MSD K.K. Approval date: 2021-12-24. Type: Partial change. Active ingredient(s): Pembrolizumab (genetical recombination).
Notes: A drug with a new indication and a new dosage for the treatment of unresectable advanced or recurrent endometrial carcinoma that has progressed after cancer chemotherapy. [Orphan drug]